PMDA approval: Tivdak for Intravenous Infusion 40 mg
Applicant: Genmab K.K. Approval date: 2025-03-27. Type: New approval. Active ingredient(s): Tisotumab vedotin (genetical recombination).
Notes: A drug with a new active ingredient indicated for the treatment of advanced or recurrent cervical cancer that has progressed after cancer chemotherapy.